PMDA approval: Rozebalamin for Injection 25 mg
Applicant: Eisai Co., Ltd. Approval date: 2024-09-24. Type: New approval. Active ingredient(s): Mecobalamin.
Notes: A drug with a new indication and a new dosage in an additional dosage form for slowing the progression of functional impairment in patients with amyotrophic lateral sclerosis (ALS). [Orphan drug]